PMDA approval: Reyvow Tablets 50 mg Reyvow Tablets 100 mg
Applicant: Eli Lilly Japan K.K. Approval date: 2022-01-20. Type: New approval. Active ingredient(s): Lasmiditan succinate.
Notes: Drugs with a new active ingredient indicated for the treatment of migraine.